PMDA approval: Imbruvica Capsules 140 mg
Applicant: Janssen Pharmaceutical K.K. Approval date: 2018-07-02. Type: Partial change. Active ingredient(s): Ibrutinib.
Notes: A drug with a new additional indication for the treatment of chronic lymphocytic leukemia (including small lymphocytic lymphoma). [Orphan drug]